PMDA approval: Augtyro Capsules 40 mg
Applicant: Bristol-Myers Squibb K.K. Approval date: 2024-09-24. Type: New approval. Active ingredient(s): Repotrectinib.
Notes: A drug with a new active ingredient indicated for the treatment of ROS1 fusion gene-positive unresectable advanced or recurrent non-small-cell lung cancer.